PMDA approval: Cibinqo Tablets 50 mg Cibinqo Tablets 100 mg Cibinqo Tablets 200 mg
Applicant: Pfizer Japan Inc. Approval date: 2021-09-27. Type: New approval. Active ingredient(s): Abrocitinib.
Notes: Drugs with a new active ingredient indicated for the treatment of atopic dermatitis in patients who have not responded sufficiently to conventional treatments.